PMDA approval: Bimzelx Syringe for S.C. Injection 160 mg Bimzelx Autoinjector for S.C. Injection 160 mg
Applicant: UCB Japan Co., Ltd. Approval date: 2024-09-24. Type: Partial change. Active ingredient(s): Bimekizumab (genetical recombination).
Notes: Drugs with a new indication and a new dosage for the treatment of hidradenitis suppurativa.